PMDA approval: Rexulti Tablets 1 mg Rexulti Tablets 2 mg
Applicant: Otsuka Pharmaceutical Co., Ltd. Approval date: 2018-01-19. Type: New approval. Active ingredient(s): Brexpiprazole.
Notes: Drugs with a new active ingredient indicated for the treatment of schizophrenia.